PMDA approval: Praluent 75 mg solution for injection in pre-filled pen Praluent 150 mg solution for injection in pre-filled pen
Applicant: Sanofi K.K. Approval date: 2018-11-21. Type: Partial change. Active ingredient(s): Alirocumab (genetical recombination).
Notes: Drugs with a new additional indication and a new dosage for the treatment of familial hypercholesterolemia and hypercholesterolemia (for use only in patients who are at higher risk of developing cardiovascular events, and in whom treatment with HMG-CoA reductase inhibitors is not suitable).